PMDA approval: Epoetin Alfa BS Injection 750 syringe [JCR] Epoetin Alfa BS Injection 1500 syringe [JCR] Epoetin Alfa BS Injection 3000 syringe [JCR] Epoetin Alfa BS Injection 750 [JCR] Epoetin Alfa BS Injection 1500 [JCR] Epoetin Alfa BS Injection 3000 [JCR]
Applicant: JCR Pharmaceuticals Co., Ltd. Approval date: 2010-01-20. Type: New approval. Active ingredient(s): Epoetin kappa (genetical recombination) [epoetin alfa biosimilar 1].
Notes: Follow-on biologics indicated for the treatment of renal anemia in patients on dialysis and anemia of prematurity.